PMDA approval: Copegus Tablets 200 mg
Applicant: Chugai Pharmaceutical Co., Ltd. Approval date: 2015-03-26. Type: Partial change. Active ingredient(s): Ribavirin.
Notes: A drug with a new additional indication and a new dosage for the improvement of viremia with the concomitant use of sofosbuvir in patients with chronic hepatitis C or compensated cirrhosis type C in serogroup 2 (genotype 2). [Expedited review]